PMDA approval: Lyxumia 300 μg solution for injection
Applicant: Sanofi K.K. Approval date: 2013-06-28. Type: New approval. Active ingredient(s): Lixisenatide.
Notes: A drug with a new active ingredient indicated for the treatment of type 2 diabetes mellitus (for use only in patients who have not responded sufficiently to either treatment (1) or (2): (1) Use of sulfonylureas (including combination with biguanides) in addition to diet and exercise therapies; (2) Use of long-acting soluble insulin or intermediate-acting insulin preparations (including combination with sulfonylureas) in addition to diet and exercise therapies.